PMDA approval: E Keppra Dry Syrup 50%
Applicant: UCB Japan Co., Ltd. Approval date: 2013-06-28. Type: New approval. Active ingredient(s): Levetiracetam.
Notes: A drug with a new additional pediatric dosage in an additional dosage form indicated for use as an adjunctive therapy with other antiepileptic drugs to treat partial seizures (including secondary generalized seizures) in patients with epilepsy who have not responded sufficiently to other antiepileptic drugs.